PMDA approval: Tavalisse Tablets 100 mg Tavalisse Tablets 150 mg
Applicant: Kissei Pharmaceutical Co., Ltd. Approval date: 2022-12-23. Type: New approval. Active ingredient(s): Fostamatinib sodium hydrate.
Notes: Drugs with a new active ingredient indicated for the treatment of chronic idiopathic thrombocytopenic purpura. [Orphan drug]